PMDA approval: Suiny Tab. 100 mg (Sanwa Kagaku Kenkyusho Co., Ltd.) Beskoa Tab. 100 mg
Applicant: Kowa Pharmaceutical Co. Ltd. Approval date: 2012-09-28. Type: New approval. Active ingredient(s): Anagliptin.
Notes: Drugs with a new active ingredient indicated for the treatment of type 2 diabetes mellitus.